PMDA approval: Bevacizumab BS Intravenous Drip Infusions 100 mg “Daiichi Sankyo” Bevacizumab BS Intravenous Drip Infusions 400 mg “Daiichi Sankyo”
Applicant: Daiichi Sankyo Company, Limited. Approval date: 2019-09-20. Type: New approval. Active ingredient(s): Bevacizumab (genetical recombination) [bevacizumab biosimilar 2].
Notes: Follow-on biologics indicated for the treatment of unresectable advanced or recurrent colon or rectal cancer.